PMDA approval: Afinitor Tablets 2.5 mg Afinitor Tablets 5 mg
Applicant: Novartis Pharma K.K. Approval date: 2014-03-17. Type: Partial change. Active ingredient(s): Everolimus.
Notes: Drugs with a new additional indication and a new dosage for the treatment of unresectable or recurrent breast cancer.